PMDA approval: Epkinly Subcutaneous Injection 4 mg Epkinly Subcutaneous Injection 48 mg
Applicant: Genmab K.K. Approval date: 2023-09-25. Type: New approval. Active ingredient(s): Epcoritamab (genetical recombination).
Notes: Drugs with a new active ingredient indicated for the treatment of relapsed or refractory large B-cell lymphoma (diffuse large B-cell lymphoma, high-grade B-cell lymphoma, and primary mediastinal large B-cell lymphoma) or relapsed or refractory follicular lymphoma.